PMDA approval: Xenpozyme for I.V. Infusion 20 mg
Applicant: Sanofi K.K. Approval date: 2022-03-28. Type: New approval. Active ingredient(s): Olipudase alfa (genetical recombination).
Notes: A drug with a new active ingredient indicated for the treatment of acid sphingomyelinase deficiency. [SAKIGAKE designation, orphan drug]